PMDA approval: Ustekinumab BS 45 mg Syringe for S.C. Injection “YD”
Applicant: Yoshindo Inc. Approval date: 2024-12-27. Type: New approval. Active ingredient(s): Ustekinumab (genetical recombination) [ustekinumab biosimilar 2].
Notes: A biosimilar indicated for the treatment of the following disease in patients who have not sufficiently responded to conventional therapies: plaque psoriasis and psoriatic arthritis